PMDA approval: Prevymis Tablets 240 mg Prevymis Intravenous Infusion 240 mg
Applicant: MSD K.K. Approval date: 2024-05-17. Type: Partial change. Active ingredient(s): Letermovir.
Notes: Drugs with a new indication for the prophylaxis of cytomegalovirus disease in organ transplantation.